PMDA approval: Avigan Tablets 200 mg
Applicant: Fujifilm Toyama Chemical Co., Ltd. Approval date: 2024-06-24. Type: Partial change. Active ingredient(s): Favipiravir.
Notes: A drug with a new indication and a new dosage for the treatment of severe fever with thrombocytopenia syndrome virus infection. [Orphan drug]